PMDA approval: Herceptin Intravenous Infusion 60 Herceptin Intravenous Infusion 150
Applicant: Chugai Pharmaceutical Co., Ltd. Approval date: 2011-11-25. Type: Partial change. Active ingredient(s): Trastuzumab (genetical recombination).
Notes: Drugs with a new additional indication and a new dosage for the treatment of breast cancer with HER2 overexpression. [Public knowledge-based application after PAFSC's preliminary assessment]